PMDA approval: Mikeluna Combination Ophthalmic Solution
Applicant: Otsuka Pharmaceutical Co., Ltd. Approval date: 2016-09-28. Type: New approval. Active ingredient(s): Carteolol hydrochloride/ Latanoprost.
Notes: A new combination drug indicated for the treatment of glaucoma and ocular hypertension.